PMDA approval: Indigocarmine Injection 20 mg “Daiichi Sankyo”
Applicant: Daiichi Sankyo Co., Ltd. Approval date: 2009-09-18. Type: Partial change. Active ingredient(s): Indigocarmine.
Notes: A drug with a new route of administration, a new indication, and a new dosage for sentinel lymph node mapping in breast cancer and malignant melanoma. [Expedited review]